PMDA approval: Rifxima Tablets 200 mg
Applicant: Aska Pharmaceutical Co., Ltd. Approval date: 2024-03-26. Type: Partial change. Active ingredient(s): Rifaximin.
Notes: A drug with a new additional pediatric dosage indicated for the improvement of hyperammonemia in patients with hepatic encephalopathy. [Orphan drug]